PMDA approval: Elneopa-NF No. 1 Injection Elneopa-NF No. 2 Injection
Applicant: Otsuka Pharmaceutical Factory Inc. Approval date: 2016-07-04. Type: New approval. Active ingredient(s): N/A for this combination drug.
Notes: Combination prescription drugs with similar formulations indicated for the supplementation of water, electrolytes, calories, amino acids, vitamins, zinc, iron, copper, manganese, and iodine in patients for whom receiving oral or enteral nutrition is impossible or insufficient and the total parenteral nutrition is the only choice.